PMDA approval: Cimzia 200 mg Syringe for S.C. Injection
Applicant: UCB Japan Co., Ltd. Approval date: 2012-12-25. Type: New approval. Active ingredient(s): Certolizumab pegol (genetical recombination).
Notes: A drug with a new active ingredient indicated for the treatment of rheumatoid arthritis (including prevention of structural joint damage) in patients who have not sufficiently responded to conventional treatments.